PMDA approval: Actemra 162 mg Syringe for SC Injection Actemra 162 mg Auto-injector for SC Injection
Applicant: Chugai Pharmaceutical Co., Ltd. Approval date: 2017-06-26. Type: Partial change. Active ingredient(s): Tocilizumab (genetical recombination).
Notes: Drugs with a new dosage indicated for the treatment of rheumatoid arthritis (including prevention of structural joint damage) in patients who have not responded sufficiently to conventional treatments.